PMDA approval: Zosyn 2.25 g for Intravenous Injection Zosyn 4.5 g for Intravenous Injection
Applicant: Taiho Pharmaceutical Co., Ltd. Approval date: 2008-07-16. Type: New approval. Active ingredient(s): Tazobactam/ piperacillin hydrate.
Notes: New combination drugs indicated for the treatment of septicemia, pneumonia, pyelonephritis, and complicated cystitis.